PMDA approval: OneDuro Patch 0.84 mg OneDuro Patch 1.7 mg OneDuro Patch 3.4 mg OneDuro Patch 5 mg OneDuro Patch 6.7 mg
Applicant: Janssen Pharmaceutical K.K. Approval date: 2013-12-20. Type: Partial change. Active ingredient(s): Fentanyl.
Notes: Drugs with a new additional indication for analgesia in moderate to severe chronic pain which cannot be managed by treatments with non -opioid analgesics and weak opioid analgesics (for use only in patients who switch from an opioid analgesic).